PMDA approval: Erelsa Tablets 50 mg
Applicant: MSD K.K. Approval date: 2016-09-28. Type: New approval. Active ingredient(s): Elbasvir.
Notes: A drug with a new active ingredient indicated for the improvement of viremia in patients with chronic hepatitis C or compensated cirrhosis type C in serogroup 1 (genotype 1). [Priority review]